PMDA approval: Cosentyx for S.C. Injection 150 mg Syringe Cosentyx for S.C. Injection 150 mg
Applicant: Novartis Pharma K.K. Approval date: 2015-12-21. Type: Partial change. Active ingredient(s): Secukinumab (genetical recombination).
Notes: Drugs with a new additional indication for the treatment of pustular psoriasis in patients who have not responded sufficiently to conventional therapies.